PMDA approval: Nesp Injection 5 μg Plastic Syringe
Applicant: Kyowa Hakko Kirin Co., Ltd. Approval date: 2013-09-20. Type: New approval. Active ingredient(s): Darbepoetin alfa (genetical recombination).
Notes: A drug with a new additional pediatric dosage in an additional dosage form. The drug is indicated for the treatment of renal anemia.